PMDA approval: Fujichenon Granular Tablets 125
Applicant: Fujimoto Pharmaceutical Corporation. Approval date: 2025-09-19. Type: New approval. Active ingredient(s): Chenodeoxycholic acid.
Notes: A drug with a new indication and a new dosage in an additional dosage form for the treatment of cerebrotendinous xanthomatosis. [Orphan drug]